PMDA approval: Magsent Injection 100 mL Magnesol for Intravenous Injection 20 mL Magsent Injection Syringe 40 mL
Applicant: Toa Pharmaceuticals Co., Ltd. Approval date: 2013-03-25. Type: Partial change. Active ingredient(s): Magnesium sulfate hydrate/glucose.
Notes: Drugs with a new additional indication and a new dosage for the prevention and treatment of eclampsia in patients with severe pregnancy-induced hypertension. [Public knowledge-based application after PAFSC's preliminary assessment]